PMDA approval: Cravit Intravenous Drip Infusion 500 mg/100 mL Cravit Intravenous Drip Infusion 500 mg/20 mL
Applicant: Daiichi Sankyo Company, Limited. Approval date: 2015-09-24. Type: Partial change. Active ingredient(s): Levofloxacin hydrate.
Notes: Drugs with new additional indications for the treatment of cystitis, pyelonephritis, secondary infection of trauma, burns or surgical wounds, and others.